PMDA approval: Valixa Dry Syrup 5000 mg
Applicant: Mitsubishi Tanabe Pharma Corporation. Approval date: 2023-03-27. Type: Partial change. Active ingredient(s): Valganciclovir hydrochloride.
Notes: A drug with a new indication and a new dosage for the treatment of symptomatic congenital cytomegalovirus infection. [Orphan drug]